PMDA approval: Effexor SR Capsules 37.5 mg Effexor SR Capsules 75 mg
Applicant: Pfizer Japan Inc. Approval date: 2015-09-28. Type: New approval. Active ingredient(s): Venlafaxine hydrochloride.
Notes: Drugs with a new active ingredient indicated for the treatment of depression.